PMDA approval: Strattera Capsule 5 mg Strattera Capsule 10 mg Strattera Capsule 25 mg Strattera Capsule 40 mg
Applicant: Eli Lilly Japan K.K. Approval date: 2012-08-24. Type: Partial change. Active ingredient(s): Atomoxetine hydrochloride.
Notes: Drugs with a new additional indication and a new dosage for the treatment of attention-deficit/hyperactivity disorder (AD/HD) in adults.